PMDA approval: Erbitux Injection 100 mg
Applicant: Merck Serono Co., Ltd. Approval date: 2012-12-21. Type: Partial change. Active ingredient(s): Cetuximab (genetical recombination).
Notes: A drug with a new additional indication for the treatment of head and neck cancer. [Priority review]